PMDA approval: Humira 40 mg for S.C. Injection Syringe 0.8 mL
Applicant: Abbott Japan Co., Ltd. Approval date: 2012-08-10. Type: Partial change. Active ingredient(s): Adalimumab (genetical recombination).
Notes: A drug with a new additional indication for the treatment of rheumatoid arthritis (including prevention of structural joint damage).